PMDA approval: Entresto Tablets 50 mg Entresto Tablets 100 mg Entresto Tablets 200 mg
Applicant: Novartis Pharma K.K. Approval date: 2024-02-09. Type: Partial change. Active ingredient(s): Sacubitril valsartan sodium hydrate.
Notes: Drugs with a new indication and a new additional pediatric dosage for the treatment of chronic heart failure.